PMDA approval: Empaveli for Subcutaneous Ingection 1080 mg
Applicant: Swedish Orphan Biovitrum Japan Co., Ltd. Approval date: 2023-03-27. Type: New approval. Active ingredient(s): Pegcetacoplan.
Notes: A drug with a new active ingredient indicated for the treatment of paroxysmal nocturnal haemoglobinuria.